PMDA approval: Proemend for Intravenous Infusion 150 mg
Applicant: Ono Pharmaceutical Co., Ltd. Approval date: 2011-09-26. Type: New approval. Active ingredient(s): Fosaprepitant meglumine.
Notes: A drug with a new active ingredient indicated for the treatment of gastrointestinal symptoms (nausea and vomiting) associated with administration of antineoplastic drugs (cisplatin, etc.) (including delayed phase).